PMDA approval: Tramcet Combination Tablets
Applicant: Janssen Pharmaceutical K.K. Approval date: 2011-04-22. Type: New approval. Active ingredient(s): Tramadol hydrochloride/ acetaminophen.
Notes: A new combination drug indicated for analgesia of chronic non-cancer pain and pain after tooth extraction which are not managed by treatments with non-opioid analgesics.